PMDA approval: Rifadin Capsules 150 mg (Daiichi Sankyo Company, Limited) Rifampicin Capsules 150 mg "Sandoz"
Applicant: Sandoz K.K. Approval date: 2011-05-20. Type: Partial change. Active ingredient(s): Rifampicin.
Notes: Drugs with a new additional indication and a new dosage for the treatment of nontuberculous mycobacteriosis including Mycobacterium avium complex (MAC) infection. [Public knowledge-based application after PAFSC's preliminary assessment]